PMDA approval: Stelues Suspension Liquid for Intramuscular Injection 600,000 Units Stelues Suspension Liquid for Intramuscular Injection 2,400,000 Units
Applicant: Pfizer Japan Inc. Approval date: 2021-09-27. Type: New approval. Active ingredient(s): Benzylpenicillin benzathine hydrate.
Notes: Drugs with a new route of administration indicated for the treatment of syphilis (excluding neurosyphilis) caused by Treponema pallidum.